PMDA approval: Vitrakvi Capsules 25 mg Vitrakvi Capsules 100 mg Vitrakvi Oral Solution 20 mg/mL
Applicant: Bayer Yakuhin, Ltd. Approval date: 2021-03-23. Type: New approval. Active ingredient(s): Larotrectinib sulfate.
Notes: Drugs with a new active ingredient indicated for the treatment of NTRK fusion gene-positive advanced or recurrent solid tumors. [Orphan drug]